PMDA approval: Entresto Tablets 50 mg Entresto Tablets 100 mg Entresto Tablets 200 mg
Applicant: Novartis Pharma K.K. Approval date: 2020-06-29. Type: New approval. Active ingredient(s): Sacubitril valsartan sodium hydrate.
Notes: Drugs with a new active ingredient indicated for the treatment of chronic heart failure (for use only in patients receiving standard treatment of chronic heart failure).